PMDA approval: Lunabell Tablets ULD
Applicant: Nobelpharma Co., Ltd. Approval date: 2013-06-28. Type: New approval. Active ingredient(s): Norethisterone/ ethinylestradiol.
Notes: A drug with a new indication and a new dosage in an additional dosage form indicated for the treatment of dysmenorrhea.